PMDA approval: Singulair Tablets 5 mg Singulair Tablets 10 (Banyu Pharmaceutical Co., Ltd.) Kipres Tablets 5 mg Kipres Tablets 10
Applicant: Kyorin Pharmaceutical Co., Ltd. Approval date: 2008-01-25. Type: New approval. Active ingredient(s): Montelukast sodium.
Notes: Drugs with a new indication and dosage and in a new dosge form for the treatment of allergic rhinitis.